PMDA approval: Paraplatin Injection 50 mg Paraplatin Injection 150 mg Paraplatin Injection 450 mg
Applicant: Bristol-Myers K.K. Approval date: 2011-11-25. Type: Partial change. Active ingredient(s): Carboplatin.
Notes: Drugs with a new additional indication and new dosages for the treatment of breast cancer. [Public knowledge-based application after PAFSC's preliminary assessment]